PMDA approval: Rituxan Intravenous Infusion 100 mg Rituxan Intravenous Infusion 500 mg
Applicant: Zenyaku Kogyo Co., Ltd. Approval date: 2024-11-22. Type: Partial change. Active ingredient(s): Rituximab (genetical recombination).
Notes: Drugs with a new additional pediatric dosage indicated for the treatment of chronic idiopathic thrombocytopenic purpura. [Public knowledge-based application after PAFSC's preliminary assessment]